PMDA approval: Faslodex Intramuscular Injection 250 mg
Applicant: AstraZeneca K.K. Approval date: 2024-03-26. Type: Partial change. Active ingredient(s): Fulvestrant.
Notes: A drug with a new dosage indicated for the treatment of breast cancer.